PMDA approval: (1) Pegintron Powder for Injection 50 μg/0.5 mL Pegintron Powder for Injection 100 μg/0.5 mL Pegintron Powder for Injection 150 μg/0.5 mL (2) Rebetol Capsules 200 mg
Applicant: MSD K.K. Approval date: 2011-12-22. Type: Partial change. Active ingredient(s): (1) Peginterferon alfa-2b (genetical recombination) (2) Ribavirin.
Notes: Drugs with a new additional indication and a new dosage for the improvement of viremia in patients with compensated cirrhosis C by concomitant use. [Priority review]